PMDA approval: Cymbalta Capsules 20 mg Cymbalta Capsules 30 mg
Applicant: Shionogi & Co., Ltd. Approval date: 2012-02-22. Type: Partial change. Active ingredient(s): Duloxetine hydrochloride.
Notes: Drugs with a new additional indication for the treatment of pain associated with diabetic neuropathy.